PMDA approval: Proemend for Intravenous Infusion 150 mg
Applicant: Ono Pharmaceutical Co., Ltd. Approval date: 2016-03-18. Type: Partial change. Active ingredient(s): Fosaprepitant meglumine.
Notes: A drug with new additional pediatric dosages indicated for the treatment of gastrointestinal symptoms (nausea and vomiting, including delayed phase) associated with administration of antineoplastic drugs (cisplatin, etc.).